PMDA approval: Leucovorin Tablets 5 mg
Applicant: Pfizer Japan Inc. Approval date: 2021-11-25. Type: Partial change. Active ingredient(s): Calcium folinate.
Notes: A drug with a new dosage indicated for the alleviation of toxicity of antifolates.